PMDA approval: Jcovden Intramuscular Injection
Applicant: Janssen Pharmaceutical K.K. Approval date: 2022-06-20. Type: New approval. Active ingredient(s): COVID-19 (SARS-CoV- 2) vaccine (recombinant adenovirus vector).
Notes: A drug with a new active ingredient indicated for the prevention of disease caused by SARS-CoV-2 infection (COVID-19). [Priority review]